PMDA approval: Volibris Tablets 2.5 mg
Applicant: GlaxoSmithKline K.K. Approval date: 2021-03-23. Type: Partial change. Active ingredient(s): Ambrisentan.
Notes: A drug with a new additional pediatric dosage indicated for the treatment of pulmonary arterial hypertension. [Orphan drug]